PMDA approval: Metopirone Capsules 250 mg
Applicant: Novartis Pharma K.K. Approval date: 2011-11-25. Type: Partial change. Active ingredient(s): Metyrapone.
Notes: A drug with a new additional indication and a new dosage for the treatment of Cushing's syndrome. [Public knowledge-based application after PAFSC's preliminary assessment]